PMDA approval: Arnuity 100 μg Ellipta 14 doses Arnuity 100 μg Ellipta 30 doses Arnuity 200 μg Ellipta 14 doses Arnuity 200 μg Ellipta 30 doses
Applicant: GlaxoSmithKline K.K. Approval date: 2017-03-30. Type: New approval. Active ingredient(s): Fluticasone furoate.
Notes: Drugs with a new dosage and other characteristics indicated for the treatment of bronchial asthma.